PMDA approval: Covgoze Intramuscular Injection
Applicant: Shionogi & Co., Ltd. Approval date: 2024-06-24. Type: New approval. Active ingredient(s): Recombinant coronavirus (SARS-CoV-2) spike protein antigen.
Notes: A drug with a new active ingredient indicated for the prevention of disease caused by SARS-CoV-2 infection (COVID-19). [Priority review]